PMDA approval: Mektovi Tablets 15 mg
Applicant: Ono Pharmaceutical Co., Ltd. Approval date: 2020-11-27. Type: Partial change. Active ingredient(s): Binimetinib.
Notes: A drug with a new indication and a new dosage for the treatment of unresectable, advanced or recurrent BRAF mutation-positive colorectal cancer that has progressed after cancer chemotherapy. [Priority review]